PMDA approval: Onetram Tablets 100 mg
Applicant: Nippon Shinyaku Co., Ltd. Approval date: 2015-03-26. Type: New approval. Active ingredient(s): Tramadol hydrochloride.
Notes: A drug in a new dosage form indicated for analgesia in patients with cancers-associated pain or chronic pain, which cannot be managed by treatments with non-opioid analgesics.